PMDA approval: Actair 100 IR Sublingual Tablets-HDM Actair 300 IR Sublingual Tablets-HDM
Applicant: Shionogi & Co., Ltd. Approval date: 2015-03-26. Type: New approval. Active ingredient(s): Dermatophagoides farinae extract bulk powder, Dermatophagoides pteronyssinus extract bulk powder.
Notes: Drugs with new active ingredients indicated for the allergen immunotherapy for house dust mite antigen-induced allergic rhinitis.